PMDA approval: Ameparomo Capsules 250 mg
Applicant: Pfizer Japan Inc. Approval date: 2012-12-25. Type: New approval. Active ingredient(s): Paromomycin sulfate.
Notes: A drug with a new active ingredient indicated for the treatment of intestinal amoebiasis.